PMDA approval: Caprelsa Tablets 100 mg
Applicant: AstraZeneca K.K. Approval date: 2015-09-28. Type: New approval. Active ingredient(s): Vandetanib.
Notes: A drug with a new active ingredient indicated for the treatment of unresectable medullary thyroid cancer. [Orphan drug]